PMDA approval: Dupixent 300 mg Syringe for S.C. Injection Dupixent 300 mg Pen for S.C. Injection
Applicant: Sanofi K.K. Approval date: 2023-06-26. Type: Partial change. Active ingredient(s): Dupilumab (genetical recombination).
Notes: Drugs with a new indication and a new dosage for the treatment of prurigo nodularis in patients who have not responded sufficiently to conventional treatments.